PMDA approval: Topiloric Tablets 20 mg Topiloric Tablets 40 mg Topiloric Tablets 60 mg (Fujiyakuhin Co., Ltd.) Uriadec Tab. 20 mg Uriadec Tab. 40 mg Uriadec Tab. 60 mg
Applicant: Sanwa Kagaku Kenkyusho Co., Ltd. Approval date: 2013-06-28. Type: New approval. Active ingredient(s): Topiroxostat.
Notes: Drugs with a new active ingredient indicated for the treatment of gout and hyperuricemia.